PMDA approval: Airwin for Subcutaneous Injection 45 mg Airwin for Subcutaneous Injection 60 mg
Applicant: MSD K.K. Approval date: 2025-06-24. Type: New approval. Active ingredient(s): Sotatercept (genetical recombination).
Notes: Drugs with a new active ingredient indicated for the treatment of pulmonary arterial hypertension. [Orphan drug]